PMDA approval: Tysabri for I.V. Infusion 300 mg
Applicant: Biogen Idec Japan Ltd. Approval date: 2014-03-24. Type: New approval. Active ingredient(s): Natalizumab (genetical recombination).
Notes: A drug with a new active ingredient indicated for the prevention of relapse and for delaying the accumulation of physical disability in multiple sclerosis. [Orphan drug]